PMDA approval: Comclo Shampoo 0.05%
Applicant: Maruho Co., Ltd. Approval date: 2021-02-24. Type: Partial change. Active ingredient(s): Clobetasol propionate.
Notes: A drug with a new indication for the treatment of eczema/dermatitis of the scalp.